PMDA approval: Rebetol Capsules 200 mg
Applicant: MSD K.K. Approval date: 2019-01-08. Type: Partial change. Active ingredient(s): Ribavirin.
Notes: A drug with a new additional indication and a new dosage, used in combination with sofosbuvir and velpatasvir, for the improvement of viremia in patients with chronic hepatitis C or compensated cirrhosis type C who have previously been treated. [Expedited review]